PMDA approval: Raiatt MIBG-I 131 Injection
Applicant: Fujifilm Toyama Chemical Co., Ltd. Approval date: 2021-09-27. Type: New approval. Active ingredient(s): 3-Iodobenzylguanidine (131I).
Notes: A drug with a new active ingredient indicated for the treatment of unresectable MIBG-positive pheochromocytoma/paraganglioma. [Orphan drug]